PMDA approval: Dobutrex Injection 100 mg Dobutrex Kit for Intravenous Infusion 200 mg Dobutrex Kit for Intravenous Infusion 600 mg (Kyowa Pharmaceutical Industry Co., Ltd.) Dobutamine Injection 100 mg [Pfizer] Dobutamine Injection 200 mg Kit [Pfizer] Dobutamine Injection 600 mg Kit [Pfizer]
Applicant: Mylan Seiyaku Ltd. Approval date: 2018-09-21. Type: Partial change. Active ingredient(s): Dobutamine hydrochloride.
Notes: Drugs with a new additional indication and a new dosage for stress echocardiography. [Public knowledge-based application after PAFSC's preliminary assessment]